PMDA approval: Rybrevant Intravenous Infusion 350 mg
Applicant: Janssen Pharmaceutical K.K. Approval date: 2025-05-19. Type: Partial change. Active ingredient(s): Amivantamab (genetical recombination).
Notes: A drug with a new dosage indicated for the treatment of unresectable advanced or recurrent epidermal growth factor receptor (EGFR ) mutation-positive non-small cell lung cancer.